PMDA approval: Lonasen Tape 20 mg Lonasen Tape 30 mg Lonasen Tape 40 mg
Applicant: Sumitomo Dainippon Pharma Co., Ltd. Approval date: 2019-06-18. Type: New approval. Active ingredient(s): Blonanserin.
Notes: Drugs with a new route of administration indicated for the treatment of schizophrenia.